PMDA approval: Gardasil Aqueous Suspension for Intramuscular Injection Syringes
Applicant: MSD K.K. Approval date: 2020-12-25. Type: Partial change. Active ingredient(s): Recombinant adsorbed 4-valent human papillomavirus virus-like particle vaccine (yeast origin).
Notes: A drug with a new indication for the prevention of the following diseases caused by infection with human papillomavirus types 6, 11, 16, and 18: • Cervical cancer (squamous cell carcinoma and adenosquamous carcinoma) and its precursor lesions (cervical intraepithelial neoplasia [CIN] 1, 2, and 3, and adenocarcinoma in situ [AIS]) • Vulvar intraepithelial neoplasia (VIN) 1, 2, and 3, and vaginal intraepithelial noeplasia (VaIN) 1, 2, and 3 • Anal cancer (squamous cell carcinoma) and its precursor lesions (anal intraepithelial neoplasia [AIN] 1, 2, and 3) • Condyloma acuminatum